PMDA approval: Tarlige Tablets 2.5 mg Tarlige Tablets 5 mg Tarlige Tablets 10 mg Tarlige Tablets 15 mg
Applicant: Daiichi Sankyo Company, Limited. Approval date: 2019-01-08. Type: New approval. Active ingredient(s): Mirogabalin besilate.
Notes: Drugs with a new active ingredient indicated for the treatment of peripheral neuropathic pain.